PMDA approval: Pomalyst Capsules 1 mg Pomalyst Capsules 2 mg Pomalyst Capsules 3 mg Pomalyst Capsules 4 mg
Applicant: Celgene K.K. Approval date: 2019-05-22. Type: Partial change. Active ingredient(s): Pomalidomide.
Notes: Drugs with a new dosage indicated for the treatment of relapsed or refractory multiple myeloma. [Orphan drug]